PMDA approval: Difolta Injection 20 mg
Applicant: Mundipharma K.K. Approval date: 2017-07-03. Type: New approval. Active ingredient(s): Pralatrexate.
Notes: A drug with a new active ingredient indicated for the treatment of relapsed or refractory peripheral T-cell lymphoma. [Orphan drug]